PMDA approval: Atozet Combination Tablets LD Atozet Combination Tablets HD
Applicant: MSD K.K. Approval date: 2017-09-27. Type: New approval. Active ingredient(s): Ezetimibe/ Atorvastatin calcium hydrate.
Notes: New combination drugs indicated for the treatment of hypercholesterolemia and familial hypercholesterolemia.